PMDA approval: Pariet Tablets 10 mg Pariet Tablets 20 mg
Applicant: Eisai Co., Ltd. Approval date: 2010-12-21. Type: Partial change. Active ingredient(s): Sodium rabeprazole.
Notes: Drugs with new dosages for the treatment of reflux esophagitis.